PMDA approval: Epogin Subcutaneous Injection Syringe 24000
Applicant: Chugai Pharmaceutical Co., Ltd. Approval date: 2010-06-18. Type: New approval. Active ingredient(s): Epoetin beta (genetical recombination).
Notes: A drug with a new dosage in a new additional dosage form indicated for autologous blood storage.